PMDA approval: Selenica-R Granules 40% Selenica-R Tab.200 mg Selenica-R Tab.400 mg
Applicant: Kowa Company, Ltd. Approval date: 2011-06-16. Type: Partial change. Active ingredient(s): Sodium valproate.
Notes: Drugs with a new additional indication and a new dosage for prevention of migraine attack.